PMDA approval: Pulmicort 100 μg Turbuhaler 112 doses Pulmicort 200 μg Turbuhaler 56 doses Pulmicort 200 μg Turbuhaler 112 doses
Applicant: AstraZeneca K.K. Approval date: 2010-07-23. Type: Partial change. Active ingredient(s): Budesonide.
Notes: Drugs with a new additional pediatric dosage indicated for the treatment of bronchial asthma.